PMDA approval: Surepost Tablets 0.25 mg Surepost Tablets 0.5 mg
Applicant: Dainippon Sumitomo Pharma Co., Ltd. Approval date: 2011-01-21. Type: New approval. Active ingredient(s): Repaglinide.
Notes: Drugs with a new active ingredient indicated for the improvement of postprandial changes of blood glucose in type 2 diabetes mellitus (for use only in patients who have not responded sufficiently to either [1] diet and exercise therapies alone or [2] alpha-glucosidase inhibitors along with diet and exercise therapies).